PMDA approval: Juxtapid Capsules 5 mg Juxtapid Capsules 10 mg Juxtapid Capsules 20 mg
Applicant: Aegerion Pharmaceuticals K.K. Approval date: 2016-09-28. Type: New approval. Active ingredient(s): Lomitapide mesilate.
Notes: Drugs with a new active ingredient indicated for the treatment of homozygous familial hypercholesterolemia. [Orphan drug]